PMDA approval: Preminent Tablets HD
Applicant: MSD K.K. Approval date: 2013-09-20. Type: New approval. Active ingredient(s): (1) Losartan potassium (2) Hydrochlorothiazide.
Notes: A drug with a new dosage in an additional dosage form. The drug is indicated for the treatment of hypertension.